PMDA approval: Hemlibra s.c. 30 mg Hemlibra s.c. 60 mg Hemlibra s.c. 90 mg Hemlibra s.c. 105 mg Hemlibra s.c. 150 mg
Applicant: Chugai Pharmaceutical Co., Ltd. Approval date: 2018-03-23. Type: New approval. Active ingredient(s): Emicizumab (genetical recombination).
Notes: Drugs with a new active ingredient indicated for the control of bleeding tendency in patients with congenital blood coagulation factor VIII deficiency with blood coagulation factor VIII inhibitors. [Orphan drug]